PMDA approval: Forteo for S.C. Injection Kit 600 μg
Applicant: Eli Lilly Japan K.K. Approval date: 2011-05-20. Type: Partial change. Active ingredient(s): Teriparatide (genetical recombination).
Notes: A drug with a new dosage with a revised treatment period from "up to 18 months" to "up to 24 months" indicated for the treatment of osteoporosis with an increased risk for fracture.